PMDA approval: Erizas Capsule for Nasal Spray 400 µg
Applicant: Nippon Shinyaku Co., Ltd. Approval date: 2009-10-16. Type: New approval. Active ingredient(s): Dexamethasone cipecilate.
Notes: A drug with a new active ingredient indicated for the treatment of allergic rhinitis.